PMDA approval: Ocnobel Tablets 150 mg Ocnobel Tablets 300 mg Ocnobel Oral Suspension 6%
Applicant: Nobelpharma Co., Ltd. Approval date: 2016-07-04. Type: New approval. Active ingredient(s): Oxcarbazepine.
Notes: Drugs with a new active ingredient indicated for the use as an adjunctive therapy with other antiepileptic drugs to treat partial seizures (including secondary generalized seizures) in patients with epilepsy who have not responded sufficiently to other antiepileptic drugs.